PMDA approval: Enjaymo for I.V. Infusion 1.1 g
Applicant: Sanofi K.K. Approval date: 2022-06-20. Type: New approval. Active ingredient(s): Sutimlimab (genetical recombination).
Notes: A drug with a new active ingredient indicated for the treatment of cold agglutinin disease. [Orphan drug]